PMDA approval: Cravit Ophthalmic Solution 1.5%
Applicant: Santen Pharmaceutical Co., Ltd. Approval date: 2010-12-21. Type: New approval. Active ingredient(s): Levofloxacin hydrate.
Notes: A drug with a new dosage of the 1.5% formulation for the treatment of blepharitis, dacryocystitis, hordeolum, conjunctivitis, meibomianitis, and keratitis (including corneal ulcer) and sterilization during the ophthalmic perioperative period.